PMDA approval: Kiklin Capsules 250 mg
Applicant: Astellas Pharma Inc. Approval date: 2012-03-30. Type: New approval. Active ingredient(s): Bixalomer.
Notes: A drug with a new active ingredient indicated for the improvement of hyperphosphatemia in patients with chronic renal failure under dialysis.